PMDA approval: Remifentanil for Intravenous Injection 2 mg "Daiichi Sankyo" Remifentanil for Intravenous Injection 5 mg "Daiichi Sankyo"
Applicant: Maruishi Pharmaceutical Co., Ltd. Approval date: 2022-08-24. Type: Partial change. Active ingredient(s): Remifentanil hydrochloride.
Notes: Drugs with a new indication and a new dosage for analgesia during artificial respiration in the intensive care setting.